PMDA approval: Calonal Fine Gran. 20% Calonal Fine Gran. 50% Calonal Tab. 200 Calonal Tab. 300 Calonal Powder (Showa Yakuhin Kako Co., Ltd.) Cocarl Dry Syr. 40% Cocarl Tab. 200 mg (Sanwa Kagaku Kenkyusho Co., Ltd.) Calsil Fine Granules 20% Calsil Tablets 200 (Taiyo Pharmaceutical Industry Co., Ltd.) Anyrume Fine Granules 20% Anyrume Tablets 200 mg Anyrume Tablets 300 mg Pyrinazin Powder (Choseido Pharmaceutical Co., Ltd.) Napa
Applicant: Mylan Seiyaku Ltd. Approval date: 2011-01-21. Type: Partial change. Active ingredient(s): Acetaminophen.
Notes: Drugs with a new additional indication for the treatment of osteoarthritis and with an expanded dosage of acetaminophen.